PMDA approval: Irbetan Tablets 50 mg Irbetan Tablets 100 mg (Shionogi & Co., Ltd.) Avapro Tablets 50 mg Avapro Tablets 100 mg
Applicant: Dainippon Sumitomo Pharma Co., Ltd. Approval date: 2008-04-16. Type: New approval. Active ingredient(s): Irbesartan.
Notes: Drugs containing a new active ingredient indicated for the treatment of hypertension.